PMDA approval: Daichirona for Intramuscular Injection
Applicant: Daiichi Sankyo Company, Limited. Approval date: 2025-03-27. Type: Partial change. Active ingredient(s): mRNA encoding the spike protein of SARS-CoV-2.
Notes: A drug with a new additional pediatric dosage indicated for the prevention of disease caused by SARS-CoV-2 infection (COVID-19).